PMDA approval: Rethio 100 mg for Intravenous Infusion
Applicant: Sumitonmo Dainippon Pharma Co., Ltd. Approval date: 2020-03-25. Type: Partial change. Active ingredient(s): Thiotepa.
Notes: A drug with a new indication and a new dosage for the conditioning treatment of malignant lymphoma in patients prior to autologous hematopoietic stem-cell transplantation.